PMDA approval: Beselna Cream 5%
Applicant: Mochida Pharmaceutical Co., Ltd. Approval date: 2007-07-31. Type: New approval. Active ingredient(s): Imiquimod.
Notes: A drug containing a new active ingredient indicated for the treatment of condyloma acuminatum (limited to the external genitals or perianal region).